PMDA approval: Ora-Bliss Gargle Gran. 11% (Showa Yakuhin Kako Co., Ltd.) Miranor Granule 11%
Applicant: Oriental Pharmaceutical and Synthetic Chemical Co., Ltd. Approval date: 2013-08-20. Type: Partial change. Active ingredient(s): Sodium fluoride.
Notes: Drugs with a new dosage indicated for the prevention of dental caries.